PMDA approval: (1) Tafinlar Dispersible Tablets for Pediatric 10 mg (2) Tafinlar Capsules 50 mg (3) Tafinlar Capsules 75 mg
Applicant: Novartis Pharma K.K. Approval date: 2024-09-24. Type: New approval. Active ingredient(s): Dabrafenib mesilate.
Notes: (1) A drug with a new indication and a new dosage in an additional dosage form for the treatment of BRAF mutation-positive low-grade glioma. (2) (3) Drugs with a new indication for the treatment of BRAF mutation-positive low-grade glioma. [Orphan drug]